PMDA approval: Rybelsus Tablets 3 mg Rybelsus Tablets 7 mg Rybelsus Tablets 14 mg
Applicant: Novo Nordisk Pharma Ltd. Approval date: 2020-06-29. Type: New approval. Active ingredient(s): Semaglutide (genetical recombination).
Notes: Drugs with a new route of administration indicated for the treatment of type 2 diabetes mellitus.